PMDA approval: Fentos Tape 1 mg Fentos Tape 2 mg Fentos Tape 4 mg Fentos Tape 6 mg Fentos Tape 8 mg
Applicant: Hisamitsu Pharmaceutical Co., Inc. Approval date: 2014-06-20. Type: Partial change. Active ingredient(s): Fentanyl citrate.
Notes: Drugs with a new indication for analgesia in moderate to severe chronic pain which cannot be managed by treatments with non-opioid analgesics and weak opioid analgesics (for use only in patients who switch from an opioid analgesic).